PMDA approval: Yervoy Injection 50 mg
Applicant: Bristol-Myers Squibb K.K. Approval date: 2020-09-25. Type: Partial change. Active ingredient(s): Ipilimumab (genetical recombination).
Notes: A drug with a new indication for the treatment of unresectable advanced or recurrent microsatellite instability-high (MSI-High) colorectal cancer that has progressed after cancer chemotherapy.